PMDA approval: Intuniv Tablets 1 mg Intuniv Tablets 3 mg
Applicant: Shionogi & Co., Ltd. Approval date: 2019-06-18. Type: Partial change. Active ingredient(s): Guanfacine hydrochloride.
Notes: Drugs with a new additional indication and a new dosage for the treatment of attention-deficit/hyperactivity disorder (AD/HD) in adults.